PMDA approval: Humira 40 mg for S.C. Injection Syringe 0.4 mL Humira 80 mg for S.C. Injection Syringe 0.8 mL Humira 40 mg for S.C. Injection in pre-filled pen 0.4 mL Humira 80 mg for S.C. Injection in pre-filled pen 0.8 mL
Applicant: AbbVie GK. Approval date: 2019-02-21. Type: Partial change. Active ingredient(s): Adalimumab (genetical recombination).
Notes: Drugs with a new additional indication and a new dosage for the treatment of hidradenitis suppurativa. [Orphan drug]